PMDA approval: Meropen for Intravenous Drip Infusion Vial 0.25 g Meropen for Intravenous Drip Infusion Vial 0.5 g Meropen for Intravenous Drip Infusion Kit 0.5 g
Applicant: Dainippon Sumitomo Pharma Co., Ltd. Approval date: 2010-01-20. Type: Partial change. Active ingredient(s): Meropenem hydrate.
Notes: Drugs with a new additional indication and a new dosage for the treatment of febrile neutropenia.